PMDA approval: Poteligeo Injection 20 mg
Applicant: Kyowa Hakko Kirin Co., Ltd. Approval date: 2012-03-30. Type: New approval. Active ingredient(s): Mogamulizumab (genetical recombination).
Notes: A drug with a new active ingredient indicated for the treatment of recurrent or relapsed CCR4-positive adult T-cell leukemia/lymphoma. [Orphan drug]